PMDA approval: Faslodex Intramuscular Injection 250 mg
Applicant: AstraZeneca K.K. Approval date: 2017-09-27. Type: Partial change. Active ingredient(s): Fulvestrant.
Notes: A drug with a revised indication and a new dosage for the treatment of breast cancer. [Expedited review]